PMDA approval: Nobelzin Capsules 25 mg Nobelzin Capsules 50 mg Nobelzin Tablets 25 mg Nobelzin Tablets 50 mg
Applicant: Nobelpharma Co., Ltd. Approval date: 2017-03-24. Type: Partial change. Active ingredient(s): Zinc acetate dihydrate.
Notes: Drugs with a new additional indication and a new dosage for the treatment of hypozincemia.